PMDA approval: Dormicum Injection 10 mg
Applicant: Astellas Pharma Inc. Approval date: 2010-03-12. Type: Partial change. Active ingredient(s): Midazolam.
Notes: A drug with a new additional pediatric dosage indicated for anesthetic premedication and for sedation during artificial respiration in patients under intensive care.